PMDA approval: Uptravi Tablets 0.2 mg Uptravi Tablets 0.4 mg
Applicant: Nippon Shinyaku Co., Ltd. Approval date: 2016-09-28. Type: New approval. Active ingredient(s): Selexipag.
Notes: Drugs with a new active ingredient indicated for the treatment of pulmonary arterial hypertension. [Orphan drug]